PMDA approval: Zithromac SR Dry Syrup 2 g for Adults
Applicant: Pfizer Japan Inc. Approval date: 2009-01-21. Type: New approval. Active ingredient(s): Azithromycin hydrate.
Notes: A drug with a new additional indication for Neisseria gonorrhoeae infections, with a new dosage in a new dosage form. Conversion from an immediate release formulation of azithromycin to a sustained release formulation.